PMDA approval: Cabpirin Combination Tablets
Applicant: Takeda Pharmaceutical Company Limited. Approval date: 2020-03-25. Type: New approval. Active ingredient(s): Aspirin/Vonoprazan fumarate.
Notes: A new combination drug indicated for inhibiting thrombus/embolization formation in the following diseases or post-operations (for use only in patients with a history of gastric ulcer or duodenal ulcer): • Angina pectoris (chronic stable angina, unstable angina), myocardial infarction, or ischemic cerebrovascular disease (transient ischemic attack [TIA], cerebral infarction) • Coronary artery bypass graft (CABG) or percutaneous transluminal coronary angioplasty (PTCA)